PMDA approval: Twotram Tab. 50 mg Twotram Tab. 100 mg Twotram Tab. 150 mg
Applicant: Nippon Zoki Pharmaceutical Co., Ltd. Approval date: 2020-09-25. Type: New approval. Active ingredient(s): Tramadol hydrochloride.
Notes: Drugs in a new dosage form indicated for analgesia in chronic pain that cannot be managed by treatment with non-opioid analgesics.